PMDA approval: Dupixent 300 mg Syringe for S.C. Injection
Applicant: Sanofi K.K. Approval date: 2018-01-19. Type: New approval. Active ingredient(s): Dupilumab (genetical recombination).
Notes: A drug with a new active ingredient indicated for the treatment of atopic dermatitis in patients who have not responded sufficiently to conventional treatments.